PMDA approval: Paxil Tablets 5 mg Paxil Tablets 10 mg Paxil Tablets 20 mg
Applicant: GlaxoSmithKline K.K. Approval date: 2013-11-22. Type: Partial change. Active ingredient(s): Paroxetine hydrochloride hydrate.
Notes: Drugs with a new additional indication and a new dosage for the treatment of posttraumatic stress disorder.